PMDA approval: Remitch Capsules 2.5 μg Remitch OD Tablets 2.5 μg (Toray Industries, Inc.) Nopicor Capsules 2.5 µg
Applicant: Toray Medical Co., Ltd. Approval date: 2017-09-22. Type: Partial change. Active ingredient(s): Nalfurafine hydrochloride.
Notes: Drugs with a revised indication for the improvement of pruritus in patients on peritoneal dialysis (for use only in patients who have not responded sufficiently to conventional treatments).